PMDA approval: Treprost 20 mg for injection Treprost 50 mg for injection Treprost 100 mg for injection Treprost 200 mg for injection
Applicant: Mochida Pharmaceutical Co., Ltd. Approval date: 2014-03-24. Type: New approval. Active ingredient(s): Treprostinil.
Notes: Drugs with a new active ingredient indicated for the treatment of pulmonary arterial hypertension (WHO functionalclassification; Class II, III and IV).